PMDA approval: Teriparatide BS Subcutaneous Injection Kit 600 μg "MOCHIDA"
Applicant: Mochida Pharmaceutical Co., Ltd. Approval date: 2019-09-20. Type: New approval. Active ingredient(s): Teriparatide (genetical recombination) [teriparatide biosimilar 1].
Notes: A follow-on biologic indicated for the treatment of osteoporosis with a high risk of fracture.